PMDA approval: Keytruda Injection 100 mg
Applicant: MSD K.K. Approval date: 2022-09-26. Type: Partial change. Active ingredient(s): Pembrolizumab (genetical recombination).
Notes: (1) A drug with a new indication for the treatment of advanced or recurrent cervical cancer. (2) A drug with a new indication and a new dosage for the preoperative and postoperative adjuvant treatment of hormone receptor (HR)-negative and human epidermal growth factor receptor 2 (HER2)-negative breast cancer with a high risk of recurrence.